PMDA approval: Busulfex Injection 60 mg
Applicant: Otsuka Pharmaceutical Co., Ltd. Approval date: 2020-03-25. Type: Partial change. Active ingredient(s): Busulfan.
Notes: A drug with a new indication and a new dosage for the conditioning treatment of malignant lymphoma in patients prior to autologous hematopoietic stem-cell transplantation. [Public knowledge-based application after PAFSC's preliminary assessment]